PMDA approval: Hunterase ICV Injection 15 mg
Applicant: Clinigen K.K. Approval date: 2021-01-22. Type: New approval. Active ingredient(s): Idursulfase beta (genetical recombination).
Notes: A drug with a new active ingredient indicated for the treatment of mucopolysaccharidosis type II. [Orphan drug]